PMDA approval: Rituxan Intravenous Infusion 100 mg Rituxan Intravenous Infusion 500 mg
Applicant: Zenyaku Kogyo Co., Ltd. Approval date: 2023-08-23. Type: Partial change. Active ingredient(s): Rituximab (genetical recombination).
Notes: Drugs with a new indication and a new dosage for the treatment of lupus nephritis in patients who have not responded sufficiently to conventional treatments. [Public knowledge-based application after preliminary assessment by the Pharmaceutical Affairs and Food Sanitation Council (PAFSC)]